PMDA approval: Imusera Capsules 0.5 mg (Mitsubishi Tanabe Pharma Corporation) Gilenya Capsules 0.5 mg
Applicant: Novartis Pharma K.K. Approval date: 2011-09-26. Type: New approval. Active ingredient(s): Fingolimod hydrochloride.
Notes: Drugs with a new active ingredient indicated for the prevention of relapse and for delaying the accumulation of physical disability in multiple sclerosis. [Orphan drug]